PMDA approval: Keytruda Injection 100 mg
Applicant: MSD K.K. Approval date: 2021-08-25. Type: Partial change. Active ingredient(s): Pembrolizumab (genetical recombination).
Notes: (1) A drug with a new indication for the treatment of unresectable advanced or recurrent microsatellite instability-high (MSI-High) colorectal cancer. (2) A drug with a new indication and a new dosage for the treatment of PD-L1-positive, hormone receptor-negative and HER2-negative inoperable or recurrent breast cancer.